PMDA approval: Pariet Tablets 5 mg Pariet Tablets 10 mg
Applicant: Eisai Co., Ltd. Approval date: 2017-09-22. Type: Partial change. Active ingredient(s): Rabeprazole sodium.
Notes: Drugs with a new dosage indicated for the treatment of reflux esophagitis.